PMDA approval: Patch Test Panel (S)
Applicant: Sato Pharmaceutical Co., Ltd. Approval date: 2014-12-26. Type: New approval. Active ingredient(s): N/A for this drug used for patch tests.
Notes: A drug indicated for use in patch tests to identify allergens in patients with allergic dermatitis.